PMDA approval: FDGscan Injectable
Applicant: Nihon Medi-Physics Co., Ltd. Approval date: 2023-08-23. Type: Partial change. Active ingredient(s): Fludeoxyglucose (18F).
Notes: A drug with a new indication for the diagnosis of malignant tumors (pleural mesothelioma, esophageal cancer, gastric cancer, gastrointestinal stromal tumor, hepatic cancer, biliary tract cancer, pancreatic cancer, bladder cancer, renal pelvis/ureter cancer, uterine cancer, ovarian cancer, bone soft tissue tumor, skin cancer [when the diagnosis of disease stage and metastasis/recurrence cannot be confirmed by other tests/imaging diagnosis]; thymic tumor, renal cancer, testicular tumor, thyroid cancer [when the diagnosis of metastasis/recurrence cannot be confirmed by other tests/imaging diagnosis]; visualization of bone lesion or extramedullary lesion in patients suspected of multiple myeloma or with multiple myeloma [when the presence of bone lesion or extramedullary lesion is suspected by other tests/imaging diagnosis]); and the visualization of inflammatory sites in patients suspected of cardiac sarcoidosis or with cardiac sarcoidosis. [Public knowledge-based application]